PMDA approval: Mohrus Tape 20 mg Mohrus Tape L 40 mg
Applicant: Hisamitsu Pharmaceutical Co., Inc. Approval date: 2011-02-23. Type: Partial change. Active ingredient(s): Ketoprofen.
Notes: Drugs with new additional indications for the treatment of myalgia and post-traumatic swelling/pain.